PMDA approval: Complavin Combination Tablets
Applicant: Sanofi K.K. Approval date: 2013-09-20. Type: New approval. Active ingredient(s): (1) Clopidogrel sulfate (2) Aspirin.
Notes: A new combination drug indicated for the treatment of ischemic heart diseases (acute coronary syndrome [unstable angina, non-ST-segment elevation myocardial infarction, ST-segment elevation myocardial infarction], stable angina, old myocardial infarction) to which percutaneous coronary intervention (PCI) is applicable.